PMDA approval: Cymbalta Capsules 20 mg Cymbalta Capsules 30 mg
Applicant: Shionogi & Co., Ltd. Approval date: 2010-01-20. Type: New approval. Active ingredient(s): Duloxetine hydrochloride.
Notes: Drugs with a new active ingredient indicated for the treatment of depression.